PMDA approval: Sonias Combination Tablets LD Sonias Combination Tablets HD
Applicant: Takeda Pharmaceutical Company Limited. Approval date: 2011-01-21. Type: New approval. Active ingredient(s): Pioglitazone hydrochloride/glimepiride.
Notes: New combination drugs indicated for the treatment of type 2 diabetes mellitus (only when a concomitant use of pioglitazone hydrochloride with glimepiride is deemed appropriate).